PMDA approval: Aloxi I.V. Injection 0.75 mg
Applicant: Taiho Pharmaceutical Co., Ltd. Approval date: 2010-01-20. Type: New approval. Active ingredient(s): Palonosetron hydrochloride.
Notes: A drug with a new active ingredient indicated for the treatment of digestive symptoms (nausea and vomiting, including delayed phase) resulting from the administration of antineoplastic agents (cisplatin, etc.).